PMDA approval: Sanglopor I.V. Infusion 2.5 g
Applicant: CSL Behring AG; designated marketing authorization holder, CSL Behring K.K. Approval date: 2010-05-13. Type: Partial change. Active ingredient(s): Freeze-dried pH4 treated human immunoglobulin.
Notes: A drug with a new dosage indicated for the treatment of hypogammaglobulinemia or agammaglobulinemia.